PMDA approval: Tazverik Tablets 200 mg
Applicant: Eisai Co., Ltd. Approval date: 2021-06-23. Type: New approval. Active ingredient(s): Tazemetostat hydrobromide.
Notes: A drug with a new active ingredient indicated for the treatment of relapsed or refractory EZH 2 gene mutation-positive follicular lymphoma (for use only if refractory or intolerant to standard therapies).